PMDA approval: Neoxy Tape 73.5 mg
Applicant: Hisamitsu Pharmaceutical Co., Inc. Approval date: 2013-03-25. Type: New approval. Active ingredient(s): Oxybutynin hydrochloride.
Notes: A drug with a new route of administration and new indications for the treatment of urinary urgency, urinary frequency, and urge urinary incontinence associated with overactive bladder.